PMDA approval: Kovaltry for iv injection 250 Kovaltry for iv injection 500 Kovaltry for iv injection 1000 Kovaltry for iv injection 2000 Kovaltry for iv injection 3000 Kovaltry for iv injection Kit 250 Kovaltry for iv injection Kit 500 Kovaltry for iv injection Kit 1000 Kovaltry for iv injection Kit 2000 Kovaltry for iv injection Kit 3000
Applicant: Bayer Yakuhin, Ltd. Approval date: 2016-03-28. Type: New approval. Active ingredient(s): Octocog beta (genetical recombination).
Notes: Drugs with a new active ingredient indicated for the control of bleeding tendency in patients with blood coagulation factor VIII deficiency.